PMDA approval: Twotram Tab. 50 mg Twotram Tab. 100 mg Twotram Tab. 150 mg
Applicant: Nippon Zoki Pharmaceutical Co., Ltd. Approval date: 2022-05-26. Type: Partial change. Active ingredient(s): Tramadol hydrochloride.
Notes: Drugs with a new indication for analgesia in various types of cancer with pain that cannot be managed by treatment with non-opioid analgesics.